PMDA approval: Accelio Intravenous Injection 1000 mg
Applicant: Terumo Corporation. Approval date: 2013-06-28. Type: New approval. Active ingredient(s): Acetaminophen.
Notes: A drug with a new route of administration indicated for the treatment of pain and pyrexia which cannnot be managed by oral preparations and suppositories.